PMDA approval: Tarlige Tablets 2.5 mg Tarlige Tablets 5 mg Tarlige Tablets 10 mg Tarlige Tablets 15 mg
Applicant: Daiichi Sankyo Company, Limited. Approval date: 2022-03-28. Type: Partial change. Active ingredient(s): Mirogabalin besilate.
Notes: Drugs with a new indication for the treatment of neuropathic pain.